PMDA approval: Itrizole Oral Solution 1%
Applicant: Janssen Pharmaceutical K.K. Approval date: 2011-09-26. Type: Partial change. Active ingredient(s): Itraconazole.
Notes: A drug with new indications for (1) the treatment of fungal infection, (2) the treatment of febrile neutropenia with suspected fungal infection, and (3) the prevention of deep mycosis in patients with hematologic malignancy or hematopoietic stem cell transplantation in whom neutropenia is anticipated.